PMDA approval: Elaprase 6 mg for Intravenous Infusion
Applicant: Genzyme Japan K.K. Approval date: 2007-10-04. Type: New approval. Active ingredient(s): Idursulfase (genetical recombination).
Notes: Drugs containing a new active ingredient indicated for the treatment of mucopolysaccharidosis II. [Orphan drug]